PMDA approval: Braftovi Capsules 50 mg Braftovi Capsules 75 mg
Applicant: Ono Pharmaceutical Co., Ltd. Approval date: 2024-05-17. Type: Partial change. Active ingredient(s): Encorafenib.
Notes: Drugs with new indications and a new dosage for the treatments of unresectable BRAF mutation-positive thyroid cancer that has progressed after cancer chemotherapy and unresectable BRAF mutation-positive anaplastic thyroid cancer.